PMDA approval: Micatrio Combination Tablets
Applicant: Nippon Boehringer Ingelheim Co., Ltd. Approval date: 2016-09-28. Type: New approval. Active ingredient(s): Telmisartan/ Amlodipine besilate/ Hydrochlorothiazide.
Notes: A new combination drug indicated for the treatment of hypertension.